PMDA approval: E-fen Buccal Tablets 50 μg E-fen Buccal Tablets 100 μg E-fen Buccal Tablets 200 μg E-fen Buccal Tablets 400 μg E-fen Buccal Tablets 600 μg E-fen Buccal Tablets 800 μg
Applicant: Teikoku Seiyaku Co., Ltd. Approval date: 2013-06-28. Type: New approval. Active ingredient(s): Fentanyl citrate.
Notes: Drugs with a new dosage in a new dosage form for the analgesia of breakthrough pain in patients with cancer receiving a potent opioid analgesic at a fixed time.